PMDA approval: Relvar 100 Ellipta 14 doses Relvar 100 Ellipta 30 doses Relvar 200 Ellipta 14 doses Relvar 200 Ellipta 30 doses
Applicant: GlaxoSmithKline K.K. Approval date: 2013-09-20. Type: New approval. Active ingredient(s): (1) Vilanterol trifenatate (2) Fluticasone furoate.
Notes: New combination drugs with a new active ingredient indicated for the treatment of bronchial asthma (when a combination treatment of an inhaled steroid and a long-acting beta-2 agonist is needed).